PMDA approval: Humira 40mg for S.C. Injection Syringe 0.8 mL
Applicant: Abbott Japan Co., Ltd. Approval date: 2010-10-27. Type: Partial change. Active ingredient(s): Adalimumab (genetical recombination).
Notes: A drug with new additional indications and a new dosage for the remission induction and maintenance therapy for moderate to severe active Crohn's disease (for use only in patients who have not sufficiently responded to conventional treatment).